PMDA approval: Imuran Tablets 50 mg (Aspen Japan K.K.) Azanin Tablets 50 mg
Applicant: Mitsubishi Tanabe Pharma Corporation. Approval date: 2019-02-21. Type: Partial change. Active ingredient(s): Azathioprine.
Notes: Drugs with a new additional indication for the treatment of autoimmune hepatitis. [Public knowledge-based application after preliminary assessment by the Pharmaceutical Affairs and Food Sanitation Council (PAFSC)]